PMDA approval: Adalimumab BS Subcutaneous Injection 20 mg Syringe 0.2 mL "CTNK" Adalimumab BS Subcutaneous Injection 40 mg Syringe 0.4 mL "CTNK" Adalimumab BS Subcutaneous Injection 80 mg Syringe 0.8 mL "CTNK" Adalimumab BS Subcutaneous Injection 40 mg Pen 0.4 mL "CTNK" Adalimumab BS Subcutaneous Injection 80 mg Pen 0.8 mL "CTNK"
Applicant: Nippon Kayaku Co., Ltd. Approval date: 2023-09-25. Type: New approval. Active ingredient(s): Adalimumab (genetical recombination) [adalimumab biosimilar 4].
Notes: Biosimilars indicated for the treatment of rheumatoid arthritis (including the prevention of structural joint damage), and the treatment of the following diseases in patients who have not sufficiently responded to conventional treatments: polyarticular-course juvenile idiopathic arthritis, plaque psoriasis, psoriatic arthritis, pustular psoriasis, ankylosing spondylitis, intestinal Behcet’ s disease, non-infectious intermediate uveitis, posterior uveitis, and panuveitis; the remission induction therapy and maintenance therapy for moderate to severe active Crohn's disease (for use only in patients who have not sufficiently responded to conventional therapies), and the treatment of moderate or severe ulcerative colitis (for use only in patients who have not sufficiently responded to conventional treatments).